PMDA approval: Lantus XR Inj. SoloStar
Applicant: Sanofi K.K. Approval date: 2015-07-03. Type: New approval. Active ingredient(s): Insulin glargine (genetical recombination).
Notes: A drug in a new dosage form indicated for the treatment of diabetes mellitus in cases where insulin therapy is indicated.